PMDA approval: Targretin Capsules 75 mg
Applicant: Minophagen Pharmaceutical Co., Ltd. Approval date: 2024-06-24. Type: Partial change. Active ingredient(s): Bexarotene.
Notes: A drug with a new indication for the treatment of adult T-cell leukemia/lymphoma with skin lesions.